PMDA approval: Cubicin IV 350 mg
Applicant: MSD K.K. Approval date: 2013-08-20. Type: Partial change. Active ingredient(s): Daptomycin.
Notes: A drug with a new dosage indicated for the treatment of sepsis, infective endocarditis, deep skin infection, secondary infection of trauma, burn and surgical wound, and secondary infection of erosion and ulcer caused by daptomycin-sensitive methicillin-resistant Staphylococcus aureus (MRSA).